PMDA approval: Fetroja for Intravenous Drip Infusion 1 g
Applicant: Shionogi & Co., Ltd. Approval date: 2023-11-30. Type: New approval. Active ingredient(s): Cefiderocol tosilate sulfate hydrate.
Notes: A drug with a new active ingredient indicated for the treatment of infections caused by Cefiderocol-sensitive Escherichia coli, Citrobacter spp, Klebsiella pneumoniae, Klebsiella spp, Enterobacter spp, Serratia marcescens, Proteus spp, Morganella morganii, Pseudomonas aeruginosa, Burkholderia spp, Stenotrophomonas maltophilia, and Acinetobacter spp (limited to bacterial strains resistant to carbapenem antimicrobial drugs). [Orphan drug]